PMDA approval: Privigen 10% I.V. Drip Infusion 5 g/50 mL Privigen 10% I.V. Drip Infusion 10 g/100 mL Privigen 10% I.V. Drip Infusion 20 g/200 mL
Applicant: CSL Behring K.K. Approval date: 2019-03-26. Type: New approval. Active ingredient(s): pH4-treated acidic normal human immunoglobulin.
Notes: Drugs with a new active ingredient indicated for the improvement of muscle weakness in chronic inflammatory demyelinating polyneuropathy and for inhibiting progression of motor disability due to chronic inflammatory demyelinating polyneuropathy (in the cases where patients show an improvement in their acute phase treatment).